PMDA approval: Pentasa Tablets 250
Applicant: Nisshin Kyorin Pharmaceutical Co., Ltd. Approval date: 2008-02-29. Type: Partial change. Active ingredient(s): Mesalazine.
Notes: A drug with a new dosage for use in pediatric patients for the treatment of ulcerative colitis (excluding severe patients) and Crohn’s disease.